PMDA approval: Zytiga Tablets 250 mg
Applicant: Janssen Pharmaceutical K.K. Approval date: 2014-07-04. Type: New approval. Active ingredient(s): Abiraterone acetate.
Notes: A drug with a new active ingredient indicated for the treatment of castration-resistant prostate cancer. [Priority review]